PMDA approval: Moviprep Combination Oral Solution
Applicant: Ajinomoto Pharmaceutical Co., Ltd. Approval date: 2012-12-25. Type: New approval. Active ingredient(s): N/A for this combination drug.
Notes: A combination prescription drug with similar formulations to be used for bowel cleansing as a preparation for colonoscopy and large intestine surgery.